PMDA approval: Unasyn-S for Intravenous Use 0.75 g Unasyn-S for Intravenous Use 1.5 g Unasyn-S Kit for Intravenous Use 1.5 g Unasyn-S Kit for Intravenous Use 3 g
Applicant: Pfizer Japan Inc. Approval date: 2012-08-10. Type: Partial change. Active ingredient(s): Sulbactam sodium/ampicillin sodium.
Notes: Drugs with new additional indications for Streptococcus Pneumoniae and Moraxella catarrhalis as applicable microorganisms. A new dosage has been added to enable high-dosage use for severe infections.